PMDA approval: Jebik V
Applicant: Research Foundation for Microbial Diseases of Osaka University. Approval date: 2009-02-23. Type: New approval. Active ingredient(s): Inactivated Japanese encephalitis virus (Beijing strain).
Notes: A drug containing a new active ingredient indicated for prophylaxis against Japanese encephalitis.